PMDA approval: Byclot Combination Intravenous Injection
Applicant: Kaketsuken [The Chemo-Sero-Therapeutic Research Institute]. Approval date: 2014-07-04. Type: New approval. Active ingredient(s): Freeze-dried activated human blood coagulation factor VII concentrate containing factor X.
Notes: A new combination drug with a new active ingredient indicated for prevention of bleeding in patients who have inhibitors against blood coagulation factor VIII or IX. [Orphan drug]